PMDA approval: Leqembi for Intravenous Infusion 200 mg Leqembi for Intravenous Infusion 500 mg
Applicant: Eisai Co., Ltd. Approval date: 2023-09-25. Type: New approval. Active ingredient(s): Lecanemab (genetical recombination).
Notes: Drugs with a new active ingredient indicated for slowing the progression of mild cognitive impairment and mild dementia due to Alzheimer’s disease. [Priority review]